PMDA approval: Patch test tapes “Nickel sulfate” 160 μg “Potassium dichromate” 19 μg “Cobalt chloride” 16 μg “Mercaptobenzothiazole” 61 μg “Formaldehyde” 150 μg “Thimerosal” 6.5 μg
Applicant: Sato Pharmaceutical Co., Ltd. Approval date: 2009-02-23. Type: New approval. Active ingredient(s): Nickel sulfate Potassium dichromate Cobalt chloride Mercaptobenzothiazole N-hydroxymethyl succinimide Thimerosal.
Notes: Drugs indicated for use in patch tests to identify allergens in patients with allergic dermatitis.